PMDA approval: (1) Xarelto Dry Syrup for Pediatric 51.7 mg (2) Xarelto Dry Syrup for Pediatric 103.4 mg (3) Xarelto Tablets 15 mg (4) Xarelto Tablets 10 mg (5) Xarelto Fine Granules 15 mg (6) Xarelto Fine Granules 10 mg (7) Xarelto OD Tablets 15 mg (8) Xarelto OD Tablets 10 mg
Applicant: Bayer Yakuhin, Ltd. Approval date: 2021-01-22. Type: New approval. Active ingredient(s): Rivaroxaban.
Notes: Drugs ([1-8]) with a new indication and a new additional pediatric dosage for the treatment and prevention of recurrent venous thromboembolism. In addition, drugs ([1, 2]) are in an additional dosage form.